PMDA approval: Alhemo Subcutaneous Injection 15 mg Alhemo Subcutaneous Injection 60 mg Alhemo Subcutaneous Injection 150 mg Alhemo Subcutaneous Injection 300 mg
Applicant: Novo Nordisk Pharma Ltd. Approval date: 2024-06-24. Type: Partial change. Active ingredient(s): Concizumab (genetical recombination).
Notes: Drugs with a new indication for the control of bleeding tendency in patients with congenital haemophilia who do not have inhibitors against blood coagulation factor VIII or IX.